PMDA approval: Gonax 80 mg for Subcutaneous Injection Gonax 120 mg for Subcutaneous Injection
Applicant: Astellas Pharma Inc. Approval date: 2012-06-29. Type: New approval. Active ingredient(s): Degarelix acetate.
Notes: Drugs with a new active ingredient indicated for the treatment of prostate cancer.